PMDA approval: Xarelto Tablets 15 mg Xarelto Tablets 10 mg
Applicant: Bayer Yakuhin, Ltd. Approval date: 2012-01-18. Type: New approval. Active ingredient(s): Rivaroxaban.
Notes: Drugs with a new active ingredient indicated for the prevention of ischemic stroke and systemic embolism in patients with nonvalvular atrial fibrillation.